PMDA approval: Protecadin Tablet 5 Protecadin Tablet 10
Applicant: Taiho Pharmaceutical Co., Ltd. Approval date: 2010-03-12. Type: Partial change. Active ingredient(s): Lafutidine.
Notes: Drugs with a new additional indication for the treatment of reflux esophagitis.